PMDA approval: Remicade for I.V. Infusion 100
Applicant: Mitsubishi Tanabe Pharma Corporation. Approval date: 2017-05-18. Type: Partial change. Active ingredient(s): Infliximab (genetical recombination).
Notes: A drug with a new dosage indicated for the treatment of Crohn’s disease. [Orphan drug]